PMDA approval: Fostoin 750mg for injection
Applicant: Nobelpharma Co., Ltd. Approval date: 2011-07-01. Type: New approval. Active ingredient(s): Fosphenytoin sodium hydrate.
Notes: A drug with a new active ingredient indicated for: 1. treatment of status epilepticus 2. prevention of seizures occurring in connection with neurosurgery, and/or consciousness disorder (head trauma, etc.) 3. temporary substitution of oral phenytoin therapy in epilepsy patients.